PMDA approval: Comirnaty Intramuscular Injection for 5 to 11 years old
Applicant: Pfizer Japan Inc. Approval date: 2022-08-30. Type: Partial change. Active ingredient(s): Tozinameran.
Notes: A drug with a new dosage indicated for the prevention of disease caused by SARS-CoV-2 infection (COVID- 19). [Drug pertaining to Special Approval for Emergency]